PMDA approval: Tagrisso Tablets 40 mg Tagrisso Tablets 80 mg
Applicant: AstraZeneca K.K. Approval date: 2016-03-28. Type: New approval. Active ingredient(s): Osimertinib mesilate.
Notes: Drugs with a new active ingredient indicated for the treatment of inoperable or recurrent epidermal growth factor receptor (EGFR) T790M mutation-positive non-small-cell lung cancer with resistance to EGFR tyrosine kinase inhibitors.